PMDA approval: Uplizna for Intravenous Infusion 100 mg
Applicant: Mitsubishi Tanabe Pharma Corporation. Approval date: 2021-03-23. Type: New approval. Active ingredient(s): Inebilizumab (genetical recombination).
Notes: A drug with a new active ingredient indicated for the prevention of relapse of neuromyelitis optica spectrum disorder (including neuromyelitis optica). [Orphan drug]